PMDA approval: Adalat-CR Tablets 10 Adalat-CR Tablets 20 Adalat-CR Tablets 40
Applicant: Bayer Yakuhin, Ltd. Approval date: 2013-06-14. Type: Partial change. Active ingredient(s): Nifedipine.
Notes: Drugs with a new dosage indicated for the treatment of hypertension.